PMDA approval: Silgard 9 Aqueous Suspension for Intramuscular Injection Syringes
Applicant: MSD K.K. Approval date: 2025-08-25. Type: Partial change. Active ingredient(s): Recombinant adsorbed 9-valent human papillomavirus virus-like particle vaccines (yeast origin).
Notes: A drug with a new indication for the prevention of the following diseases caused by infection with human papillomavirus types 6, 11, 16, 18, 31, 33, 45, 52, and 58: ・Anal cancer (squamous cell carcinoma) and its precursor lesions (anal intraepithelial neoplasia [AIN] 1, 2, and 3)